PMDA approval: Geninax Tablets 200 mg
Applicant: Toyama Chemical Co., Ltd. Approval date: 2007-07-31. Type: New approval. Active ingredient(s): Garenoxacin mesilate.
Notes: A drug containing a new active ingredient indicated for the treatment of laryngopharyngitis, tonsillitis (including peritonsillitis and peritonsillar abscess), acute bronchitis, pneumonia, infections secondary to chronic respiratory disease, otitis media, and sinusitis.